PMDA approval: Vpriv for Intravenous Injection 400 U
Applicant: Shire Japan KK. Approval date: 2014-07-04. Type: New approval. Active ingredient(s): Velaglucerase alfa (genetical recombination).
Notes: A drug with a new active ingredient indicated for the improvement of various symptoms of Gaucher disease (anemia, thrombocytopenia, hepatosplenomegaly, and bone disease). [Orphan drug]